PMDA approval: Remicade for I.V. Infusion 100
Applicant: Mitsubishi Tanabe Pharma Corporation. Approval date: 2010-06-18. Type: Partial change. Active ingredient(s): Infliximab (genetical recombination).
Notes: A drug with a new additional indication and a new dosage for the treatment of moderate to severe ulcerative colitis (for use only in patients who have not sufficiently responded to conventional treatments).